PMDA approval: MenQuadfi intramuscular injection
Applicant: Sanofi K.K. Approval date: 2022-09-26. Type: New approval. Active ingredient(s): Neisseria meningitidis polysaccharides conjugated to tetanus toxoid (serogroups A, C, W, and Y).
Notes: A drug with a new active ingredient indicated for the prophylaxis of invasive meningococcal disease caused by Neisseria meningitidis (serogroups A, C, W and Y).